PMDA approval: Rexulti Tablets 1 mg Rexulti Tablets 2 mg Rexulti OD Tablets 0.5 mg Rexulti OD Tablets 1 mg Rexulti OD Tablets 2 mg
Applicant: Otsuka Pharmaceutical Co., Ltd. Approval date: 2024-09-24. Type: Partial change. Active ingredient(s): Brexpiprazole.
Notes: Drugs with new indications and a new dosage for the treatment of excessive motor activity or verbal/physical aggression attributable to rapid changes in mood and/or irritability, outburst associated with Alzheimer's dementia.